PMDA approval: Trastuzumab BS for Intravenous Infusion 60 mg [Pfizer] Trastuzumab BS for Intravenous Infusion 150 mg [Pfizer]
Applicant: Pfizer Japan Inc. Approval date: 2018-09-21. Type: New approval. Active ingredient(s): Trastuzumab (genetical recombination) [trastuzumab biosimilar 3].
Notes: Follow-on biologics indicated for the treatment of breast cancer overexpressing HER2 and unresectable advanced or recurrent gastric cancer overexpressing HER2.